PMDA approval: Lyxumia 300 μg solution for injection
Applicant: Sanofi K.K. Approval date: 2016-12-19. Type: Partial change. Active ingredient(s): Lixisenatide.
Notes: A drug with a revised indication for the treatment of type 2 diabetes mellitus.